PMDA approval: Daichirona for Intramuscular Injection
Applicant: Daiichi Sankyo Company, Limited. Approval date: 2023-11-28. Type: Partial change. Active ingredient(s): Ufrenmeran, MAFB- 7256a.
Notes: A drug with a new indication, a new dosage, and other characteristics for the prevention of disease caused by SARS-CoV-2 infection (COVID-19).